PMDA approval: Pulmicort Respules 0.25 mg Pulmicort Respules 0.5 mg
Applicant: AstraZeneca K.K. Approval date: 2010-11-16. Type: Partial change. Active ingredient(s): Budesonide.
Notes: Drugs with new additional adult and pediatric (5 years of age and older) dosages for the treatment of bronchial asthma.